PMDA approval: Akalux IV Infusion 250 mg
Applicant: Rakuten Medical Japan K.K. Approval date: 2020-09-25. Type: New approval. Active ingredient(s): Cetuximab sarotalocan sodium (genetical recombination).
Notes: A drug with a new active ingredient indicated for the treatment of unresectable locally advanced or recurrent head and neck cancer. [SAKIGAKE designation, Conditional early approval]